PMDA approval: Pemazyre Tablets 4.5 mg
Applicant: Incyte Biosciences Japan G.K. Approval date: 2023-03-27. Type: Partial change. Active ingredient(s): Pemigatinib.
Notes: A drug with a new indication and a new dosage for the treatment of FGFR1 fusion gene-positive myeloid/lymphoid neoplasms. [Orphan drug]